PMDA approval: Allelock Granule 0.5%
Applicant: Kyowa Hakko Kirin Co., Ltd. Approval date: 2011-07-01. Type: New approval. Active ingredient(s): Olopatadine hydrochloride.
Notes: A drug with an additional dosage form of granules and with a new additional pediatric dosage indicated for the treatment of allergic rhinitis, urticaria, and itching associated with skin disease (eczema/dermatitis and pruritus cutaneous).